PMDA approval: kenketu Glovenin-I for I.V. Injection 2500 mg kenketu Glovenin-I for I.V. Injection 500 mg kenketu Glovenin-I for I.V. Injection 5000 mg
Applicant: Nihon Pharmaceutical Co., Ltd. Approval date: 2014-07-04. Type: Partial change. Active ingredient(s): Freeze-dried polyethylene glycol treated human normal immunoglobulin.
Notes: Drugs with new additional indications for the treatment of Stevens-Johnson syndrome and toxic epidermal necrolysis (for use when steroid drugs are not sufficiently effective ). [Orphan drug]